PMDA approval: Taltz 80mg Syringe for SC Injection Taltz 80mg Auto-Injector for SC Injection
Applicant: Eli Lilly Japan K.K. Approval date: 2016-07-04. Type: New approval. Active ingredient(s): Ixekizumab (genetical recombination).
Notes: Drugs with a new active ingredient indicated for the treatment of plaque psoriasis, psoriatic arthritis, pustular psoriasis, and erythrodermic psoriasis in patients who have not responded sufficiently to conventional therapies.